PMDA approval: Lunesta Tablets 1 mg Lunesta Tablets 2 mg Lunesta Tablets 3 mg
Applicant: Eisai Co., Ltd. Approval date: 2012-01-18. Type: New approval. Active ingredient(s): Eszopiclone.
Notes: Drugs with a new active ingredient indicated for the treatment of insomnia.